PMDA approval: Doptelet Tablets 20 mg
Applicant: Swedish Orphan Biovitrum Japan Co., Ltd. Approval date: 2023-03-27. Type: New approval. Active ingredient(s): Avatrombopag maleate.
Notes: A drug with a new active ingredient indicated for the improvement of thrombocytopenia in patients with chronic liver disease for whom an elective invasive procedure is planned.